PMDA approval: Dovobet Ointment
Applicant: Leo Pharma K.K. Approval date: 2014-07-04. Type: New approval. Active ingredient(s): (1) Calcipotriol hydrate/ (2) Betamethasone dipropionate.
Notes: A new combination drug indicated for the treatment of psoriasis vulgaris.